PMDA approval: Emend Capsules 80 mg Emend Capsules 125 mg Emend Capsules Set
Applicant: Ono Pharmaceutical Co., Ltd. Approval date: 2009-10-16. Type: New approval. Active ingredient(s): Aprepitant.
Notes: Drugs with a new active ingredient indicated for the treatment of digestive symptoms (nausea and vomiting, including delayed phase) resulting from the administration of antineoplastic agents (cisplatin, etc.).